PMDA approval: Kytril Injection 1 mg Kytril Injection 3 mg Kytril Intravenous Bag 3 mg/50 mL Kytril Intravenous Bag 3 mg/100 mL
Applicant: Taiyo Pharma Co., Ltd. Approval date: 2022-02-25. Type: Partial change. Active ingredient(s): Granisetron hydrochloride.
Notes: Drugs with a new indication and a new dosage for the treatment of digestive symptoms (nausea and vomiting) after surgery. [Public knowledge-based application after PAFSC's preliminary assessment]